PMDA approval: Salagen Tablets 5 mg
Applicant: Kissei Pharmaceutical Co., Ltd. Approval date: 2007-10-19. Type: Partial change. Active ingredient(s): Pilocarpine hydrochloride.
Notes: A drug with a new indication for the treatment of oral dryness in patients with Sjögren’s syndrome.